PMDA approval: Synflorix Aqueous Suspension for Intramuscular Injection
Applicant: Japan Vaccine Co., Ltd. Approval date: 2015-03-26. Type: New approval. Active ingredient(s): Pneumococcal 10-valent conjugate vaccine adsorbed (Non-Typeable Haemophilus influenzae (NTHi) protein D, diphtheria or tetanus toxoid conjugates).
Notes: A drug with a new active ingredient indicated for the prophylaxis of pneumonia and pneumococcal invasive disease. (serotypes 1, 4, 5, 6B, 7F, 9V, 14, 18C, 19F, and 23F)